PMDA approval: Exjade Dispersible Tablets 125 mg Exjade Dispersible Tablets 500 mg
Applicant: Novartis Pharma K.K. Approval date: 2008-04-16. Type: New approval. Active ingredient(s): Deferasirox.
Notes: Drugs with a new active ingredient indicated for the treatment of chronic iron overload due to blood transfusions (in patients for whom injection of iron chelating agents is inappropriate). [Priority review]